PMDA approval: Yervoy Injection 20 mg Yervoy Injection 50 mg
Applicant: Bristol-Myers Squibb K.K. Approval date: 2022-05-26. Type: Partial change. Active ingredient(s): Ipilimumab (genetical recombination).
Notes: Drugs with a new indication and a new dosage for the treatment of unresectable advanced or recurrent esophageal cancer. [Priority review]